PMDA approval: Recarbrio Combination for Intravenous Drip Infusion
Applicant: MSD K.K. Approval date: 2021-06-23. Type: New approval. Active ingredient(s): Relebactam hydrate/Imipenem hydrate/Cilastatin sodium.
Notes: A new combination drug with a new active ingredient indicated for the treatment of infections caused by Recarbrio-sensitive Escherichia coli, Citrobacter spp., Klebsiella spp., Enterococcus spp., Serratia spp., Pseudomonas aeruginosa, and Acinetobacter spp. (limited to the bacterial strains resistant to carbapenem antimicrobial drugs) [Orphan drug]